PMDA approval: Pombiliti for I.V. Infusion 105 mg
Applicant: Amicus Therapeutics, Inc. Approval date: 2025-06-24. Type: New approval. Active ingredient(s): Cipaglucosidase alfa (genetical recombination).
Notes: A drug with a new active ingredient indicated for use as a combination therapy with miglustat for the treatment of late-onset Pompe disease (LOPD). [Orphan drug]